PMDA approval: Endoxan for Injection 100 mg Endoxan for Injection 500 mg
Applicant: Shionogi & Co., Ltd. Approval date: 2019-03-26. Type: Partial change. Active ingredient(s): Cyclophosphamide hydrate.
Notes: Drugs with other characteristics indicated for the treatment prior to tumor-specific T-cell infusion therapy. [Expedited review]